PMDA approval: Symbicort Turbuhaler 30 doses Symbicort Turbuhaler 60 doses
Applicant: AstraZeneca K.K. Approval date: 2009-10-16. Type: New approval. Active ingredient(s): Budesonide/formoterol fumarate hydrate.
Notes: New combination drugs indicated for the treatment of bronchial asthma (when a combination treatment of an inhaled steroid and a long-acting beta-2 agonist is needed).